PMDA approval: NovoEight i.v.injection 250 NovoEight i.v.injection 500 NovoEight i.v.injection 1000 NovoEight i.v.injection 1500 NovoEight i.v.injection 2000 NovoEight i.v.injection 3000
Applicant: Novo Nordisk Pharma Ltd. Approval date: 2014-01-17. Type: New approval. Active ingredient(s): Turoctocog alpha (genetical recombination).
Notes: Drugs with a new active ingredient indicated for inhibition of bleeding tendency in patients with blood coagulation factor VIII deficiency.